PMDA approval: Hernicore 1.25 Units for Intradiscal Inj.
Applicant: Seikagaku Corporation. Approval date: 2018-03-23. Type: New approval. Active ingredient(s): Condoliase.
Notes: A drug with a new active ingredient indicated for the treatment of lumbar disc herniation with subligamentous extrusion that has not adequately responded to conservative treatment.